PMDA approval: Vaxneuvance Aqueous Suspension Syringes
Applicant: MSD K.K. Approval date: 2023-06-26. Type: Partial change. Active ingredient(s): Pneumococcal polysaccharides of serotypes 1, 3, 4, 5, 6A, 6B, 7F, 9V, 14, 18C, 19A, 19F, 22F, 23F, and 33F conjugated to CRM197 carrier protein.
Notes: A drug with a new indication and a new additional pediatric dosage for the prophylaxis of pneumococcal disease (serotypes 1, 3, 4, 5, 6A, 6B, 7F, 9V, 14, 18C, 19A, 19F, 22F, 23F, and 33F) in individuals who are considered to be at a high risk of pneumococcal disease and invasive pneumococcal disease (serotypes 1, 3, 4, 5, 6A, 6B, 7F, 9V, 14, 18C, 19A, 19F, 22F, 23F and 33F) in children.